PMDA approval: Actair 100 IR Sublingual Tablets-HDM Actair 300 IR Sublingual Tablets-HDM
Applicant: Shionogi & Co., Ltd. Approval date: 2018-02-16. Type: Partial change. Active ingredient(s): Dermatophagoides farinae extract bulk powder, Dermatophagoides pteronyssinus extract bulk powder.
Notes: Drugs with a new additional pediatric dosage for children aged under 12 years. These drugs comprise allergen immunotherapy for the treatment of house dust mite antigen-induced allergic rhinitis.